PMDA approval: Provocholine Powder for Inhalation Solution 100 mg (Sanwa Kagaku Kenkyusho Co., Ltd.) Kenbran Powder for Inhalation Solution 100 mg
Applicant: Santen Pharmaceutical Co., Ltd. Approval date: 2016-01-22. Type: New approval. Active ingredient(s): Methacholine chloride.
Notes: Drugs with a new active ingredient indicated for tests in the diagnosis of bronchial airway hyperresponsiveness.